PMDA approval: Velcade Injection 3 mg
Applicant: Janssen Pharmaceutical K.K. Approval date: 2018-03-23. Type: Partial change. Active ingredient(s): Bortezomib.
Notes: A drug with a new additional indication and a new dosage indicated for the treatment of Waldenströ m's macroglobulinemia and lymphoplasmacytic lymphoma. [Public knowledge-based application after PAFSC's preliminary assessment]